PMDA approval: Velcade Injection 3 mg
Applicant: Janssen Pharmaceutical K.K. Approval date: 2015-06-26. Type: Partial change. Active ingredient(s): Bortezomib.
Notes: A drug with a new additional indication and a new dosage for the treatment of patients with mantle cell lymphoma. [Orphan drug]